PMDA approval: Opdivo Intravenous Infusion 20 mg Opdivo Intravenous Infusion 100 mg Opdivo Intravenous Infusion 120 mg Opdivo Intravenous Infusion 240 mg
Applicant: Ono Pharmaceutical Co., Ltd. Approval date: 2024-02-09. Type: Partial change. Active ingredient(s): Nivolumab (genetical recombination).
Notes: Drugs with a new indication and a new dosage for the treatment of unresectable advanced or recurrent epithelial skin malignancies. [Orphan drug]